PMDA approval: Habekacin Injection 25 mg Habekacin Injection 75 mg Habekacin Injection 100 mg Habekacin Injection 200 mg
Applicant: Meiji Seika Kaisha, Ltd. Approval date: 2008-02-29. Type: Partial change. Active ingredient(s): Arbekacin sulfate.
Notes: Drugs with a new dosage and in a new dosage form indicated for the treatment of septicemia and pneumonia caused by arbekacin-sensitive methicillin-resistant staphylococcus aureus (MRSA).